PMDA approval: Sirturo Tablets 100 mg
Applicant: Janssen Pharmaceutical K.K. Approval date: 2018-01-19. Type: New approval. Active ingredient(s): Bedaquiline fumarate.
Notes: A drug with a new active ingredient indicated for the treatment of pulmonary multidrug-resistant tuberculosis. [Orphan drug]